PMDA approval: Forxiga 5 mg Tablets Forxiga 10 mg Tablets
Applicant: AstraZeneca K.K. Approval date: 2021-08-25. Type: Partial change. Active ingredient(s): Dapagliflozin propylene glycolate hydrate.
Notes: Drugs with a new indication for the treatment of chronic kidney disease. [Priority review]